PMDA approval: Fentos Tape 0.5 mg Fentos Tape 1 mg Fentos Tape 2 mg Fentos Tape 4 mg Fentos Tape 6 mg Fentos Tape 8 mg
Applicant: Hisamitsu Pharmaceutical Co., Inc. Approval date: 2018-07-02. Type: New approval. Active ingredient(s): Fentanyl citrate.
Notes: Drugs with a new dosage and in additional dosage form indicated for: (1) analgesia in various types of cancer with moderate to severe pain that cannot be managed with non-opioid analgesics or weak opioid analgesics (for use only in patients who switch from other opioid analgesics); or (2) analgesia in moderate to severe chronic pain (for use only in patients who switch from other opioid analgesics).